PMDA approval: Afinitor Tablets 5 mg Afinitor Tablets 2.5 mg
Applicant: Novartis Pharma K.K. Approval date: 2016-08-26. Type: Partial change. Active ingredient(s): Everolimus.
Notes: Drugs with a revised indication from "pancreatic neuroendocrine tumor" to "neuroendocrine tumor."